PMDA approval: Bridion Intravenous 200 mg Bridion Intravenous 500 mg
Applicant: Schering-Plough K.K. Approval date: 2010-01-20. Type: New approval. Active ingredient(s): Sugammadex sodium.
Notes: Drugs with a new active ingredient indicated for the recovery from neuromuscular blockade induced by rocuronium bromide or vecuronium bromide.